PMDA approval: Soliris for Intravenous Infusion 300 mg
Applicant: Alexion Pharma G.K. Approval date: 2013-09-13. Type: Partial change. Active ingredient(s): Eculizumab (genetical recombination).
Notes: A drug with a new additional indication and a new dosage for the treatment of patients with atypical hemolytic uremic syndrome (aHUS) to inhibit complement-mediated thrombotic microangiopathy.